PMDA approval: Prograf Capsules 0.5 mg Prograf Capsules 1 mg
Applicant: Astellas Pharma Inc. Approval date: 2013-06-14. Type: Partial change. Active ingredient(s): Tacrolimus hydrate.
Notes: Drugs with a new additional indication and new dosage for the treatment of interstitial pneumonia associated with polymyositis/dermatomyositis. [Orphan drug]